PMDA approval: Cyanokit Injection Set
Applicant: Merck Ltd., Japan. Approval date: 2007-09-28. Type: New approval. Active ingredient(s): Hydroxocobalamin.
Notes: Drugs with a new indication and dosage for the treatment of cyanogen and cyanide poisoning.